PMDA approval: Sarclisa 100 mg I.V. Infusion Sarclisa 500 mg I.V. Infusion
Applicant: Sanofi K.K. Approval date: 2025-02-20. Type: Partial change. Active ingredient(s): Isatuximab (genetical recombination).
Notes: Drugs with a new indication and a new dosage for the treatment of multiple myeloma.